PMDA approval: Prolia Subcutaneous Injection 60 mg Syringe
Applicant: Daiichi Sankyo Company, Limited. Approval date: 2017-07-03. Type: Partial change. Active ingredient(s): Denosumab (genetical recombination).
Notes: A drug with a new additional indication and a new dosage indicated for inhibiting progression of bone erosion associated with rheumatoid arthritis.